PMDA approval: Telavic 250 mg Tablet
Applicant: Mitsubishi Tanabe Pharma Corporation. Approval date: 2014-09-19. Type: Partial change. Active ingredient(s): Telaprevir.
Notes: A drug with a new additional indication for the improvement of viremia in serogroup 2 (genotype III [2a] or IV [2b]) chronic hepatitis C in patients who did not respond to, or in whom the symptom relapsed with, interferon monotherapy or interferon-ribavirin combination therapy. [Priority review]